PMDA approval: Pulmozyme Inhalation Solution 2.5 mg
Applicant: Chugai Pharmaceutical Co., Ltd. Approval date: 2012-03-30. Type: New approval. Active ingredient(s): Dornase alfa (genetical recombination).
Notes: A drug with a new active ingredient indicated for the improvement of lung function in patients with cystic fibrosis. [Orphan drug]